PMDA approval: Cylocide Injection 20 mg Cylocide Injection 40 mg Cylocide Injection 60 mg Cylocide Injection 100 mg Cylocide Injection 200 mg
Applicant: Nippon Shinyaku Co., Ltd. Approval date: 2023-05-25. Type: Partial change. Active ingredient(s): Cytarabine.
Notes: Drugs with a new dosage indicated for the treatment of acute leukemia. [Public knowledge-based application, Expedited review]